PMDA approval: Daklinza Tablets 60 mg (Bristol-Myers K.K.) Sunvepra Capsules 100 mg
Applicant: Bristol-Myers K.K. Approval date: 2015-03-20. Type: Partial change. Active ingredient(s): Daclatasvir hydrochloride Asunaprevir.
Notes: Drugs with a revised indication for the improvement of viremia in patients with chronic hepatitis C or compensated cirrhosis type C in serogroup 1 (genotype 1). [Priority review]